PMDA approval: Levemir Penfill Levemir FlexPen Levemir InnoLet
Applicant: Novo Nordisk Pharma Ltd. Approval date: 2011-09-16. Type: New approval. Active ingredient(s): Insulin detemir (genetical recombination).
Notes: Drugs with a new active ingredient indicated for the treatment of diabetes mellitus in cases where insulin therapy is indicated. This application applies only to the change in manufacturing method of the drug substance, and thus formulation, manufacturing method, indications and dosage and administration of the product are the same as the approved product.